PMDA approval: Cortiment Tablets 9 mg
Applicant: Ferring Pharmaceuticals Co., Ltd. Approval date: 2023-06-26. Type: New approval. Active ingredient(s): Budesonide.
Notes: A drug with a new route of administration indicated for the treatment of active ulcerative colitis (excluding severe cases).